PMDA approval: Parsabiv Intravenous Injection for Dialysis 2.5 mg Parsabiv Intravenous Injection for Dialysis 5 mg Parsabiv Intravenous Injection for Dialysis 10 mg
Applicant: Ono Pharmaceutical Co., Ltd. Approval date: 2016-12-19. Type: New approval. Active ingredient(s): Etelcalcetide hydrochloride.
Notes: Drugs with a new active ingredient indicated for the treatment of secondary hyperparathyroidism in patients on hemodialysis.